PMDA approval: Narusus Tablets 2 mg Narusus Tablets 6 mg Narusus Tablets 12 mg Narusus Tablets 24 mg Narurapid Tablets 1 mg Narurapid Tablets 2 mg Narurapid Tablets 4 mg
Applicant: Daiichi Sankyo Propharma Co., Ltd. Approval date: 2017-03-30. Type: New approval. Active ingredient(s): Hydromorphone hydrochloride.
Notes: Drugs with a new active ingredient indicated for management of moderate to severe pain in various types of cancer.